PMDA approval: Opdivo Intravenous Infusion 20 mg Opdivo Intravenous Infusion 100 mg
Applicant: Ono Pharmaceutical Co., Ltd. Approval date: 2016-02-29. Type: Partial change. Active ingredient(s): Nivolumab (genetical recombination).
Notes: Drugs with a new dosage indicated for the treatment of unresectable melanoma. [Orphan drug]